PMDA approval: Melbin Tablets 250 mg (Dainippon Sumitomo Pharma Co., Ltd.) Glycoran Tablets 250 mg
Applicant: Nippon Shinyaku Co., Ltd. Approval date: 2009-05-20. Type: Partial change. Active ingredient(s): Metformin hydrochloride.
Notes: Drugs with a new indication and a new dosage for the treatment of type 2 diabetes mellitus in patients who have not responded sufficiently to either (1) diet and exercise therapies alone or (2) sulfonylurea along with diet and exercise therapies.